PMDA approval: Endoxan for Injection 100 mg Endoxan for Injection 500 mg
Applicant: Shionogi & Co., Ltd. Approval date: 2022-11-24. Type: Partial change. Active ingredient(s): Cyclophosphamide hydrate.
Notes: Drugs with a new dosage indicated for the treatment of breast cancer (preoperative or postoperative chemotherapy in patients with operable breast cancer). [Public knowledge-based application]